PMDA approval: Ezharmia Tablets 50 mg Ezharmia Tablets 100 mg
Applicant: Daiichi Sankyo Company, Limited. Approval date: 2024-06-24. Type: Partial change. Active ingredient(s): Valemetostat tosilate.
Notes: Drugs with a new indication for the treatment of relapsed or refractory peripheral T-cell lymphoma. [SAKIGAKE review]